PMDA approval: Asacol Tablets 400 mg
Applicant: Zeria Pharmaceutical Co., Ltd. Approval date: 2017-05-18. Type: Partial change. Active ingredient(s): Mesalazine.
Notes: A drug with a new dosage indicated for the treatment of ulcerative colitis (excluding severe cases).